PMDA approval: Lunabell Tablets
Applicant: Nobelpharma Co., Ltd. Approval date: 2010-12-21. Type: Partial change. Active ingredient(s): Norethisterone/ ethinylestradiol.
Notes: Drugs with a new additional indication for the treatment of functional dysmenorrhoea.